PMDA approval: Kindaly 4E Kindaly 4D Kindaly AF-4P
Applicant: Fuso Pharmaceutical Industries, Ltd. Approval date: 2010-12-21. Type: New approval. Active ingredient(s): N/A for this combination drug.
Notes: Combination prescription drugs with similar formulations to be used as the perfusate of dialysis-based artificial kidneys in chronic renal failure (use in patients whose blood sugar level is not controlled well with sugarless dialysate and when other bicarbonate dialysate is not sufficiently effective to improve hyperkalemia and hypermagnesemia, or when hypercalcemia may occur).